PMDA approval: Alhemo Subcutaneous Injection 15 mg Alhemo Subcutaneous Injection 60 mg Alhemo Subcutaneous Injection 150 mg Alhemo Subcutaneous Injection 300 mg
Applicant: Novo Nordisk Pharma Ltd. Approval date: 2023-09-25. Type: New approval. Active ingredient(s): Concizumab (genetical recombination).
Notes: Drugs with a new active ingredient indicated for the control of bleeding tendency in patients with congenital haemophilia who have inhibitors against blood coagulation factor VIII or IX. [Orphan drug]